PMDA approval: Ngenla Inj. 24 mg Pens Ngenla Inj. 60 mg Pens
Applicant: Pfizer Japan Inc. Approval date: 2022-01-20. Type: New approval. Active ingredient(s): Somatrogon (genetical recombination).
Notes: Drugs with a new active ingredient indicated for the treatment of short stature due to growth hormone deficiency prior to epiphyseal closure.